PMDA approval: Metoana Combination Tablets LD Metoana Combination Tablets HD
Applicant: Sanwa Kagaku Kenkyusho Co., Ltd. Approval date: 2018-09-21. Type: New approval. Active ingredient(s): Anagliptin/Metformin hydrochloride.
Notes: New combination drugs indicated for the treatment of type 2 diabetes mellitus (only when a concomitant use of anagliptin with metformin hydrochloride is deemed appropriate).